PMDA approval: Kindaly AF-5 Kindaly AF-5P Kindaly 5E
Applicant: Fuso Pharmaceutical Industries, Ltd. Approval date: 2020-06-29. Type: New approval. Active ingredient(s): N/A for this combination drug.
Notes: Combination prescription drugs with similar formulations to be used as perfusion fluids for hemodialysis in patients with chronic renal failure (use in any of the following cases: 1. when the patient’s blood sugar level is not controlled well with sugarless dialysate, 2. when other bicarbonate dialysate is not sufficiently effective to improve hyperkalemia, 3. when other bicarbonate dialysate is not sufficiently effective to improve hypermagnesemia, 4. when other bicarbonate dialysate may cause hypercalcemia).